PMDA approval: Upasita IV Injection 25 µg Syringe for Dialysis Upasita IV Injection 50 µg Syringe for Dialysis Upasita IV Injection 100 µg Syringe for Dialysis Upasita IV Injection 150 µg Syringe for Dialysis Upasita IV Injection 200 µg Syringe for Dialysis Upasita IV Injection 250 µg Syringe for Dialysis Upasita IV Injection 300 µg Syringe for Dialysis
Applicant: Sanwa Kagaku Kenkyusho Co., Ltd. Approval date: 2021-06-23. Type: New approval. Active ingredient(s): Upacicalcet sodium hydrate.
Notes: Drugs with a new active ingredient indicated for the treatment of secondary hyperparathyroidism in patients on hemodialysis.